PMDA approval: Co-Dio Combination Tablets MD Co-Dio Combination Tablets EX
Applicant: Novartis Pharma K.K. Approval date: 2009-01-21. Type: New approval. Active ingredient(s): Valsartan/ hydrochlorothiazide.
Notes: New combination drugs indicated for the treatment of hypertension.